PMDA approval: Symbicort Turbuhaler 30 doses Symbicort Turbuhaler 60 doses
Applicant: AstraZeneca K.K. Approval date: 2012-06-22. Type: Partial change. Active ingredient(s): Budesonide/formoterol fumarate hydrate.
Notes: Drugs with a new dosage for inhalation as needed in addition to periodic inhalation as a maintenance therapy. These drugs are indicated for the treatment of bronchial asthma (when a combination treatment of an inhaled steroid and a long-acting beta-2 agonist is needed).